PMDA approval: Inisync Combination Tablets
Applicant: Takeda Pharmaceutical Company Limited. Approval date: 2016-09-28. Type: New approval. Active ingredient(s): Alogliptin benzoate/ Metformin hydrochloride.
Notes: A new combination drug indicated for the treatment of type 2 diabetes mellitus (only when a concomitant use of alogliptin benzoate with metformin hydrochloride is deemed appropriate).